PMDA approval: Olumiant Tablets 4 mg Olumiant Tablets 2 mg
Applicant: Eli Lilly Japan K.K. Approval date: 2017-07-03. Type: New approval. Active ingredient(s): Baricitinib.
Notes: Drugs with a new active ingredient indicated for the treatment of rheumatoid arthritis (including prevention of structural joint damage) in patients who have not responded sufficiently to conventional treatments.